PMDA approval: Xeloda Tablets 300
Applicant: Chugai Pharmaceutical Co., Ltd. Approval date: 2016-08-26. Type: Partial change. Active ingredient(s): Capecitabine.
Notes: A drug with a new additional indication and a new dosage for adjuvant chemotherapy for rectal cancer. [Public knowledge-based application after PAFSC's preliminary assessment]